PMDA approval: Vyvgart for Intravenous Infusion 400 mg
Applicant: Argenx Japan K.K. Approval date: 2024-03-26. Type: Partial change. Active ingredient(s): Efgartigimod alfa (genetical recombination).
Notes: A drug with a new indication and a new dosage for the treatment of chronic idiopathic thrombocytopenic purpura. [Orphan drug]